PMDA approval: Strensiq Subcutaneous Injection 12 mg/0.3 mL Strensiq Subcutaneous Injection 18 mg/0.45 mL Strensiq Subcutaneous Injection 28 mg/0.7 mL Strensiq Subcutaneous Injection 40 mg/1 mL Strensiq Subcutaneous Injection 80 mg/0.8 mL
Applicant: Alexion Pharma G.K. Approval date: 2015-07-03. Type: New approval. Active ingredient(s): Asfotase alfa (genetical recombination).
Notes: Drugs with a new active ingredient indicated for the treatment of hypophosphatasia. [Orphan drug]